PMDA approval: Depromel Tablets 25 Depromel Tablets 50 Depromel Tablets 75 (Meiji Seika Pharma Co., Ltd.) Luvox Tablets 25 Luvox Tablets 50 Luvox Tablets 75
Applicant: AbbVie GK. Approval date: 2017-07-03. Type: Partial change. Active ingredient(s): Fluvoxamine maleate.
Notes: Drugs with a new additional pediatric dosage indicated for the treatment of obsessive-compulsive disorder.